PMDA approval: Neoral Oral Solution 10% Neoral 10 mg Capsules Neoral 25 mg Capsules Neoral 50 mg Capsules
Applicant: Novartis Pharma K.K. Approval date: 2017-08-25. Type: Partial change. Active ingredient(s): Ciclosporin.
Notes: Drugs with a revised indication and a new dosage for the treatment of aplastic anemia which is not severe. [Expedited review]